PMDA approval: Rinvoq Tablets 7.5 mg Rinvoq Tablets 15 mg
Applicant: AbbVie GK. Approval date: 2023-02-24. Type: Partial change. Active ingredient(s): Upadacitinib hydrate.
Notes: Drugs with a new indication and a new dosage for the treatment of non-radiographic axial spondyloarthritis in patients who have not responded sufficiently to conventional treatments.